PMDA approval: Tykerb Tablets 250 mg
Applicant: GlaxoSmithKline K.K. Approval date: 2009-04-22. Type: New approval. Active ingredient(s): Lapatinib tosilate hydrate.
Notes: A drug with a new active ingredient indicated for the treatment of inoperable or recurrent breast cancer with HER2 overexpression. [Priority review]